PMDA approval: Abrysvo Intramuscular Injection
Applicant: Pfizer Japan Inc. Approval date: 2024-03-26. Type: Partial change. Active ingredient(s): Respiratory syncytial virus-A prefusion F protein Respiratory syncytial virus-B prefusion F protein.
Notes: A drug with a new indication and a new dosage for the prevention of disease caused by respiratory syncytial virus infection in individuals 60 years of age and older.